PMDA approval: Plavix 25 mg Tablets Plavix 75 mg Tablets
Applicant: Sanofi-Aventis K.K. Approval date: 2012-08-24. Type: Partial change. Active ingredient(s): Clopidogrel sulfate.
Notes: Drugs with a new additional indication for the treatment of the following ischemic heart disease for which percutaneous coronary intervention (PCI) is applied: ST elevation myocardial infarction.